PMDA approval: Bilanoa Tablet 20 mg
Applicant: Taiho Pharmaceutical Co. Ltd. Approval date: 2016-09-28. Type: New approval. Active ingredient(s): Bilastine.
Notes: A drug with a new active ingredient indicated for the treatment of allergic rhinitis, urticaria, and itching associated with skin diseases (eczema/dermatitis, cutaneous pruritus).